PMDA approval: Imbruvica Capsules 140 mg
Applicant: Janssen Pharmaceutical K.K. Approval date: 2021-09-27. Type: Partial change. Active ingredient(s): Ibrutinib.
Notes: A drug with a new indication and a new dosage for the treatment of chronic graft-versus-host disease after hematopoietic stem cell transplantation (for use when steroid drugs are not sufficiently effective). [Orphan drug]